PMDA approval: Treakisym Injection 25 mg Treakisym Injection 100 mg
Applicant: SymBio Pharmaceuticals Limited. Approval date: 2019-03-26. Type: Partial change. Active ingredient(s): Bendamustine hydrochloride.
Notes: Drugs with other characteristics indicated for the treatment prior to tumor-specific T-cell infusion therapy. [Expedited review]